PMDA approval: Metoject Subcutaneous Injection 7.5 mg Syringe 0.15 mL Metoject Subcutaneous Injection 10 mg Syringe 0.20 mL Metoject Subcutaneous Injection 12.5 mg Syringe 0.25 mL Metoject Subcutaneous Injection 15 mg Syringe 0.30 mL
Applicant: nippon medac Co., Ltd. Approval date: 2022-09-26. Type: New approval. Active ingredient(s): Methotrexate.
Notes: Drugs with a new indication and a new dosage in an additional dosage form for the treatment of rheumatoid arthritis.